PMDA approval: Reblozyl for S.C. Injection 25 mg Reblozyl for S.C. Injection 75 mg
Applicant: Bristol-Myers Squibb K.K. Approval date: 2024-01-18. Type: New approval. Active ingredient(s): Luspatercept (genetical recombination).
Notes: Drugs with a new active ingredient indicated for the treatment of anemia associated with myelodysplastic syndrome. [Orphan drug]